PMDA approval: Adoair 250 Diskus
Applicant: GlaxoSmithKline K.K. Approval date: 2009-01-21. Type: Partial change. Active ingredient(s): Salmeterol xinafoate/fluticasone propionate.
Notes: A drug with a new additional indication and a new dosage for the alleviation of various symptoms associated with chronic obstructive pulmonary disease (chronic bronchitis and emphysema) (for use when concomitant use of inhaled corticosteroid and long-acting β2-agonist is required).